PMDA approval: Evusheld Intramuscular Injection Set
Applicant: AstraZeneca K.K. Approval date: 2022-08-30. Type: New approval. Active ingredient(s): Tixagevimab (genetical recombination), Cilgavimab (genetical recombination).
Notes: A drug with new active ingredients indicated for the treatment and suppression of development of disease caused by SARS-CoV-2 infection (COVID-19). [Drug pertaining to Special Approval for Emergency]